PMDA approval: Sujanu Combination Tablets
Applicant: MSD K.K. Approval date: 2018-03-23. Type: New approval. Active ingredient(s): Sitagliptin phosphate hydrate/Ipragliflozin L-proline.
Notes: A new combination drug indicated for the treatment of type 2 diabetes mellitus (only when a concomitant use of sitagliptin phosphate hydrate with ipragliflozin L-proline is deemed appropriate).